PMDA approval: E Keppra for I.V. Infusion 500 mg
Applicant: UCB Japan Co., Ltd. Approval date: 2014-07-04. Type: New approval. Active ingredient(s): Levetiracetam.
Notes: A drug with a new route of administration indicated for use as an adjunctive therapy with other antiepileptic drugs to treat partial seizure (including secondary generalized seizure) in patients with epilepsy who have not responded sufficiently to other antiepileptic drugs. It is used as an alternative therapy for levetiracetam oral formulation in patients who are temporarily unable to be administered orally.